PMDA approval: Halaven Injection 1 mg
Applicant: Eisai Co., Ltd. Approval date: 2011-04-22. Type: New approval. Active ingredient(s): Eribulin mesylate.
Notes: A drug with a new active ingredient indicated for the treatment of inoperable or recurrent breast cancer. [Priority review]